PMDA approval: Actos Tablets 15 Actos Tablets 30
Applicant: Takeda Pharmaceutical Company Limited. Approval date: 2009-03-24. Type: Partial change. Active ingredient(s): Pioglitazone hydrochloride.
Notes: Drugs with a new indication and a new dosage for the treatment of Type 2 diabetes mellitus in patients who have not benefitted sufficiently from taking insulin preparations in conjunction with dietary and exercise regimens in whom insulin resistance is suspected.